PMDA approval: Vimizim Intravenous Infusion 5 mg
Applicant: BioMarin Pharmaceutical Japan K.K. Approval date: 2014-12-26. Type: New approval. Active ingredient(s): Elosulfase alfa (genetical recombination).
Notes: A drug with a new active ingredient indicated for the treatment of mucopolysaccharidosis type IVA. [Orphan drug]